PMDA approval: Adempas Tablets 0.5 mg Adempas Tablets 1.0 mg Adempas Tablets 2.5 mg
Applicant: Bayer Yakuhin, Ltd. Approval date: 2015-02-20. Type: Partial change. Active ingredient(s): Riociguat.
Notes: Drugs with a new additional indication for the treatment of pulmonary arterial hypertension.